PMDA approval: Lumigan Ophthalmic Solution 0.03%
Applicant: Senju Pharmaceutical Co., Ltd. Approval date: 2009-07-07. Type: New approval. Active ingredient(s): Bimatoprost.
Notes: A drug with a new active ingredient indicated for the treatment of glaucoma and ocular hypertension.